PMDA approval: Praluent 75 mg solution for injection in pre-filled pen Praluent 150 mg solution for injection in pre-filled pen Praluent 75 mg solution for injection in pre-filled syringe Praluent 150 mg solution for injection in pre-filled syringe
Applicant: Sanofi K.K. Approval date: 2016-07-04. Type: New approval. Active ingredient(s): Alirocumab (genetical recombination).
Notes: Drugs with a new active ingredient indicated for the treatment of familial hypercholesteremia and hypercholesterolemia (for use only in patients who are at higher risk of developing cardiovascular event and have not responded sufficiently to HMG-CoA reductase inhibitors).